PMDA approval: Haiyitan Tablets 50 mg
Applicant: Haihe Biopharma K. K. Approval date: 2024-06-24. Type: New approval. Active ingredient(s): Gumarontinib hydrate.
Notes: A drug with a new active ingredient indicated for the treatment of unresectable advanced or recurrent MET exon 14 (METex14) skipping mutation-positive non-small cell lung cancer.